PMDA approval: Seibule Tablets 25 mg Seibule Tablets 50 mg Seibule Tablets 75 mg
Applicant: Sanwa Kagaku Kenkyusho Co., Ltd. Approval date: 2009-11-06. Type: Partial change. Active ingredient(s): Miglitol.
Notes: Drugs with a new additional indication for improvement of postprandial hyperglycemia in patients with diabetes mellitus (for use only in patients who have not responded sufficiently to treatment with biguanides in conjunction with dietary and exercise regimens).